PMDA approval: Onpattro for Intravenous Infusion 2 mg/mL
Applicant: Alnylam Japan K.K. Approval date: 2019-06-18. Type: New approval. Active ingredient(s): Patisiran sodium.
Notes: A drug with a new active ingredient indicated for the treatment of transthyretin familial amyloid polyneuropathy. [Orphan drug]